PMDA approval: Tamiflu Dry Syrup 3% Tamiflu Capsule 75
Applicant: Chugai Pharmaceutical Co., Ltd. Approval date: 2009-12-18. Type: Partial change. Active ingredient(s): Oseltamivir phosphate.
Notes: Drugs with a new additional indication and a new dosage for prophylaxis of influenza A or B virus infections.